PMDA approval: Orencia for I.V. Infusion 250 mg Orencia SC 125 mg Syringe 1 mL Orencia SC 125 mg Autoinjector 1 mL
Applicant: Bristol-Myers Squibb K.K. Approval date: 2020-02-21. Type: Partial change. Active ingredient(s): Abatacept (genetical recombination).
Notes: Drugs with a new additional indication for the treatment of rheumatoid arthritis (including the prevention of structural joint damage) in patients who have not responded sufficiently to conventional treatments.